PMDA approval: Revolade Tablets 12.5 mg Revolade Tablets 25 mg
Applicant: Novartis Pharma K.K. Approval date: 2024-11-22. Type: Partial change. Active ingredient(s): Eltrombopag olamine.
Notes: Drugs with a new additional pediatric dosage indicated for the treatment of chronic idiopathic thrombocytopenic purpura. [Public knowledge-based application after preliminary assessment by the Pharmaceutical Affairs and Food Sanitation Council (PAFSC)]